PMDA approval: (1) Dupixent 200 mg Syringe for S.C. Injection (2) Dupixent 300 mg Syringe for S.C. Injection (3) Dupixent 300 mg Pen for S.C. Injection
Applicant: Sanofi K.K. Approval date: 2023-09-25. Type: New approval. Active ingredient(s): Dupilumab (genetical recombination).
Notes: (1) A drug with a new additional pediatric dosage in an additional dosage form and (2), (3) drugs with a new additional pediatric dosage, indicated for the treatment of atopic dermatitis in patients who have not sufficiently responded to conventional treatments.